PMDA approval: Lafenta Tape 1.38 mg Lafenta Tape 2.75 mg Lafenta Tape 5.5 mg Lafenta Tape 8.25 mg Lafenta Tape 11 mg
Applicant: Nippon Zoki Pharmaceutical Co., Ltd. Approval date: 2019-03-26. Type: New approval. Active ingredient(s): Fentanyl.
Notes: Drugs in a new dosage form indicated for analgesia in various types of cancer with moderate to severe pain that cannot be managed with non-opioid analgesics or weak opioid analgesics (for use only in patients who switch from other opioid analgesics).